PMDA approval: Rybrevant Intravenous Infusion 350 mg
Applicant: Janssen Pharmaceutical K.K. Approval date: 2024-09-24. Type: New approval. Active ingredient(s): Amivantamab (genetical recombination).
Notes: A drug with a new active ingredient indicated for the treatment of unresectable advanced or recurrent epidermal growth factor receptor (EGFR) gene exon 20 insertion mutation-positive non-small cell lung cancer.